PMDA approval: Nisseki Polyglobin-N 5% I.V. 2.5 g/50 mL
Applicant: Japanese Red Cross Society. Approval date: 2010-05-13. Type: Partial change. Active ingredient(s): pH4-treated acidic human normal immunoglobulin.
Notes: A drug with a new dosage indicated for the treatment of hypogammaglobulinemia or agammaglobulinemia.